PMDA approval: Imfinzi Injection 120 mg Imfinzi Injection 500 mg
Applicant: AstraZeneca K.K. Approval date: 2020-08-21. Type: Partial change. Active ingredient(s): Durvalumab (genetical recombination).
Notes: Drugs with a new indication and a new dosage for the treatment of extensive-stage small-cell lung cancer.